PMDA approval: Oldamin for Injection 1 g
Applicant: Fuji Chemical Industries Co., Ltd. Approval date: 2024-12-27. Type: Partial change. Active ingredient(s): Monoethanolamine oleate.
Notes: A drug with a new indication and a new dosage for the use in sclerotherapy of venous malformation.